PMDA approval: Trazenta Tablets 5 mg
Applicant: Nippon Boehringer Ingelheim Co., Ltd. Approval date: 2011-07-01. Type: New approval. Active ingredient(s): Linagliptin.
Notes: A drug with a new active ingredient indicated for the treatment of type 2 diabetes mellitus (for use only in patients who have not responded sufficiently to diet and exercise therapies alone).